PMDA approval: Alecensa Capsules 150mg
Applicant: Chugai Pharmaceutical Co., Ltd. Approval date: 2024-08-28. Type: Partial change. Active ingredient(s): Alectinib hydrochloride.
Notes: A drug with a new indication, a new dosage, and other characteristics for the postoperative adjuvant treatment for anaplastic lymphoma kinase (ALK ) fusion gene-positive non-small cell lung cancer. [Orphan drug]